PMDA approval: Maintate Tablets 2.5 mg Maintate Tablets 5 mg
Applicant: Mitsubishi Tanabe Pharma Corporation. Approval date: 2013-06-14. Type: Partial change. Active ingredient(s): Bisoprolol fumarate.
Notes: Drugs with a new additional indication and a new dosage for the treatment of tachycardiac atrial fibrillation.